PMDA approval: Niflec
Applicant: Ajinomoto Co., Inc. Approval date: 2009-04-22. Type: Partial change. Active ingredient(s): N/A for this combination drug.
Notes: A drug with a new additional indication and a new dosage for cleansing of gastrointestinal tract for pretreatment of barium enema X-ray examination. Gasmotin Tablets 2.5 mg Gasmotin Tablets 5 mg Gasmotin Powder (Dainippon Sumitomo Pharma Co., Ltd.) Change Change Change Mosapride citrate hydrate Drugs with a new additional indication and a new dosage for adjunctive treatment to pretreatment with orally gastrointestinal lavage solution for barium enema X-ray examination.